PMDA approval: Canaglu Tablets 100 mg
Applicant: Mitsubishi Tanabe Pharma Corporation. Approval date: 2022-06-20. Type: Partial change. Active ingredient(s): Canagliflozin hydrate.
Notes: A drug with a new indication for the treatment of chronic kidney disease associated with type 2 diabetes mellitus (excluding patients who have end-stage renal failure or are undergoing dialysis).